PMDA approval: Vyalev Combination Subcutaneous infusion
Applicant: AbbVie GK. Approval date: 2022-12-23. Type: New approval. Active ingredient(s): Foslevodopa, Foscarbidopa hydrate.
Notes: A new combination drug with new active ingredients indicated for the improvement of wearing-off phenomenon in the symptoms of Parkinson's disease in patients who have not responded sufficiently to conventional levodopa-containing drug therapies.